PMDA approval: Cyramza Injection 100 mg Cyramza Injection 500 mg
Applicant: Eli Lilly Japan K.K. Approval date: 2016-06-20. Type: Partial change. Active ingredient(s): Ramucirumab (genetical recombination).
Notes: Drugs with a new additional indication and a new dosage for the treatment of unresectable advanced/relapsed non-small cell lung cancer.